PMDA approval: Suglat Tablets 25 mg Suglat Tablets 50 mg
Applicant: Astellas Pharma Inc. Approval date: 2014-01-17. Type: New approval. Active ingredient(s): Ipragliflozin L-proline.
Notes: Drugs with a new active ingredient indicated for the treatment of type 2 diabetes mellitus.